PMDA approval: Lynspad for Intravenous Infusion 1000 mg
Applicant: Grifols Therapeutics LLC. Approval date: 2021-01-22. Type: New approval. Active ingredient(s): Lyophilized human alpha1-proteinase inhibitor concentrate.
Notes: A drug with a new active ingredient indicated for the augmentation and maintenance therapy of severe alpha 1-antitrypsin deficiency. [Orphan drug]